PMDA approval: TS-1 Capsule 20 TS-1 Capsule 25
Applicant: Taiho Pharmaceutical Co., Ltd. Approval date: 2007-08-23. Type: Partial change.
Notes: A combination drug of Tegafur, Gimeracil, and Oteracil potassium Drugs with a new indication for the treatment of biliary cancer in addition to gastric cancer, colorectal cancer, head and neck cancer, non-small cell lung cancer, inoperable or relapsed breast cancer, and pancreatic cancer.